PMDA approval: Cell Culture-derived Influenza Vaccine H5N1 "Baxter" (Baxter Limited) Cell Culture-derived Influenza Vaccine H5N1 "Takeda" 5 mL
Applicant: Takeda Pharmaceutical Company Limited. Approval date: 2013-06-18. Type: New approval. Active ingredient(s): Cell culture-derived influenza vaccine (H5N1).
Notes: Drugs with a new active ingredient indicated for the prevention of pandemic influenza (H5N1). [Orphan drug]